PMDA approval: Xofluza Tablets 20 mg Xofluza Granules 2%
Applicant: Shionogi & Co., Ltd. Approval date: 2020-11-27. Type: Partial change. Active ingredient(s): Baloxavir marboxil.
Notes: Drugs with a new indication and a new dosage for the prophylaxis of influenza A or B virus infection.